PMDA approval: Uritos Tablets 0.1 mg (Kyorin Pharmaceutical Co., Ltd.) Staybla Tablets 0.1 mg
Applicant: Ono Pharmaceutical Co., Ltd. Approval date: 2009-12-18. Type: Partial change. Active ingredient(s): Imidafenacin.
Notes: Drugs with a new dosage indicated for the treatment of urgency of urination, pollakiuria, and urge urinary incontinence associated with overactive bladder.